PMDA approval: Abilify Tablets 3 mg Abilify Tablets 6 mg Abilify Tablets 12 mg Abilify OD Tablets 3 mg Abilify OD Tablets 6 mg Abilify OD Tablets 12 mg Abilify Powder 1% Abilify Oral Solution 0.1%
Applicant: Otsuka Pharmaceutical Co., Ltd. Approval date: 2013-06-14. Type: Partial change. Active ingredient(s): Aripiprazole.
Notes: Drugs with a new additional indication and a new dosage for the treatment of depression (for use only in patients who have an inadequate response to antidepressant therapy).